PMDA approval: Goofice Tablets 5 mg
Applicant: EA Pharma Co., Ltd. Approval date: 2018-01-19. Type: New approval. Active ingredient(s): Elobixibat hydrate.
Notes: A drug with a new active ingredient indicated for the treatment of chronic constipation (excluding constipation due to organic diseases).